PMDA approval: LenaDex Tablets 4 mg
Applicant: Celgene K.K. Approval date: 2010-06-18. Type: New approval. Active ingredient(s): Dexamethasone.
Notes: A drug with a new additional indication and a new dosage for the treatment of multiple myeloma. [Expedited review]